PMDA approval: Lipidil Tablets 53.3 mg Lipidil Tablets 80 mg (Aska Pharmaceutical Co., Ltd.) Tricor Tablets 53.3 mg Tricor Tablets 80 mg
Applicant: Abbott Japan Co., Ltd. Approval date: 2011-03-30. Type: New approval. Active ingredient(s): Fenofibrate.
Notes: Drugs in new dosage forms and with new dosages indicated for the treatment of hyperlipemia (including familial hyperlipemia).